PMDA approval: Jardiance Tablets 25 mg Jardiance Tablets 10 mg
Applicant: Nippon Boehringer Ingelheim Co., Ltd. Approval date: 2014-12-26. Type: New approval. Active ingredient(s): Empagliflozin.
Notes: Drugs with a new active ingredient indicated for the treatment of type 2 diabetes mellitus.